PMDA approval: Prevymis Tablets 240 mg Prevymis Intravenous Infusion 240 mg
Applicant: MSD K.K. Approval date: 2018-03-23. Type: New approval. Active ingredient(s): Letermovir.
Notes: Drugs with a new active ingredient indicated for the prophylaxis of cytomegalovirus disease in allogeneic hematopoietic stem cell transplant recipients. [Orphan drug]